PMDA approval: Livmarli Oral Solution 10 mg/mL
Applicant: Takeda Pharmaceutical Company Limited. Approval date: 2025-03-27. Type: New approval. Active ingredient(s): Maralixibat chloride.
Notes: A drug with a new active ingredient indicated for the treatment of cholestatic pruritus in patients with Alagille syndrome and progressive familial intrahepatic cholestasis. [Orphan drug]